PMDA approval: Mekinist Tablets 0.5 mg Mekinist Tablets 2 mg
Applicant: Novartis Pharma K.K. Approval date: 2018-07-02. Type: Partial change. Active ingredient(s): Trametinib dimethyl sulfoxide.
Notes: Drugs with a new additional indication and a new dosage indicated for the treatment of BRAF mutation-positive melanoma. [Orphan drug]